PMDA approval: Nesp Injection 10 μg Plastic Syringe Nesp Injection 15 μg Plastic Syringe Nesp Injection 20 μg Plastic Syringe Nesp Injection 30 μg Plastic Syringe Nesp Injection 40 μg Plastic Syringe Nesp Injection 60 μg Plastic Syringe Nesp Injection 120 μg Plastic Syringe Nesp Injection 180 μg Plastic Syringe
Applicant: Kyowa Hakko Kirin Co., Ltd. Approval date: 2013-09-13. Type: Partial change. Active ingredient(s): Darbepoetin alfa (genetical recombination).
Notes: Drugs with a new additional pediatric dosage. These drugs are indicated for the treatment of renal anemia.